PMDA approval: Xeloda Tablet 300
Applicant: Chugai Pharmaceutical Co., Ltd. Approval date: 2009-09-18. Type: Partial change. Active ingredient(s): Capecitabine.
Notes: A drug with a new additional indication and a new additional dosage and administration for combination therapy with other anticancer drugs (XELOX + BV regimen) for advanced or recurrent colorectal cancer not suited for curative resection.